PMDA approval: Alabel Oral 1.5 g (Nobelpharma Co., Ltd.) Alaglio Internal Medicine 1.5 g
Applicant: SBI Pharmaceuticals Co., Ltd. Approval date: 2013-03-25. Type: New approval. Active ingredient(s): Aminolevulinic acid hydrochloride.
Notes: Drugs with a new active ingredient indicated for the visualization of tumor tissues during tumorectomy for malignant glioma. [Orphan drug]